PMDA approval: Septocaine Combination Injection Cartridge
Applicant: GC Showayakuhin Corporation. Approval date: 2024-09-24. Type: New approval. Active ingredient(s): Articaine hydrochloride/ Adrenaline bitartrate.
Notes: A new combination drug with a new active ingredient indicated for infiltration anesthesia or nerve block in the fields of dentistry and oral surgery.